PMDA approval: Yervoy Injection 50 mg
Applicant: Bristol-Myers Squibb K.K. Approval date: 2020-11-27. Type: Partial change. Active ingredient(s): Ipilimumab (genetical recombination).
Notes: A drug with a new indication and a new dosage for the treatment of unresectable advanced or recurrent non-small cell lung cancer.